PMDA approval: Comirnaty Intramuscular Injection for 5 to 11 years old
Applicant: Pfizer Japan Inc. Approval date: 2022-01-21. Type: New approval. Active ingredient(s): Tozinameran.
Notes: A drug with a new pediatric dosage and a drug in an additional dosage form indicated for the prevention of disease caused by SARS-CoV-2 infection (COVID-19). [Drug pertaining to Special Approval for Emergency]